PMDA approval: NovoSeven for Injection 1.2 mg NovoSeven for Injection 4.8 mg
Applicant: Novo Nordisk Pharma Ltd. Approval date: 2010-03-12. Type: Partial change. Active ingredient(s): Eptacog alfa (activated) (genetical recombination).
Notes: Drugs with a new additional indication and a new dosage for inhibition of bleeding tendency in patients with congenital factor VII deficiency. 77 NovoSeven HI for Intravenous Injection 1 mg NovoSeven HI for Intravenous Injection 2 mg NovoSeven HI for Intravenous Injection 5 mg (Novo Nordisk Pharma Ltd.) Change Change Change